PMDA approval: Baktar Combination Tablets Baktar Combination Granules (Shionogi & Co., Ltd.) Bactramin Combination Tablet Bactramin Combination Granule
Applicant: Chugai Pharmaceutical Co., Ltd. Approval date: 2012-08-10. Type: Partial change. Active ingredient(s): Sulfamethoxazole/ trimethoprim.
Notes: Drugs with a new additional indication and a new dosage for the treatment and prevention of Pneumocystis pneumonia. [Public knowledge-based application after PAFSC's preliminary assessment]